PMDA approval: Breztri Aerosphere 56 Inhalations Breztri Aerosphere 120 Inhalations
Applicant: AstraZeneca K.K. Approval date: 2019-06-18. Type: New approval. Active ingredient(s): Budesonide /Glycopyrronium bromide/Formoterol fumarate hydrate.
Notes: New combination drugs with a new active ingredient indicated for the relief of symptoms in patients with chronic obstructive pulmonary disease (chronic bronchitis, emphysema) (who require a combination therapy with an inhaled corticosteroid, a long-acting inhaled anticholinergic agent and a long-acting beta-2 agonist).